PMDA approval: Vasolan Tablets 40 mg
Applicant: Eizai Co., Ltd. Approval date: 2008-02-29. Type: Partial change. Active ingredient(s): Verapamil hydrochloride.
Notes: A drug with a new indication for the treatment of tachyarrhythmia (atrial fibrillation and flutter, and paroxysmal supraventricular tachycardia).